PMDA approval: Genotropin TC Inj. 5.3 mg Genotropin TC Inj. 12 mg Genotropin GoQuick Inj. 5.3 mg Genotropin GoQuick Inj. 12 mg
Applicant: Pfizer Japan Inc. Approval date: 2023-12-22. Type: Partial change. Active ingredient(s): Somatropin (genetical recombination).
Notes: Drugs with a new indication and a new dosage for the improvement of body composition in patients with Prader-Willi syndrome. [Orphan drug]